PMDA approval: Dormicum Injection 10 mg
Applicant: Astellas Pharma Inc. Approval date: 2013-12-20. Type: Partial change. Active ingredient(s): Midazolam.
Notes: A drug with a new additional indication and a new dosage for sedation during surgeries and other procedures in the field of dentistry and oral surgery.